PMDA approval: Differin Gel 0.1%
Applicant: Galderma S.A. Approval date: 2008-07-16. Type: New approval. Active ingredient(s): Adapalene.
Notes: A drug containing a new active ingredient indicated for the treatment of acne vulgaris.